PMDA approval: Ofev Capsules 100 mg Ofev Capsules 150 mg
Applicant: Nippon Boehringer Ingelheim Co., Ltd. Approval date: 2019-12-20. Type: Partial change. Active ingredient(s): Nintedanib ethanesulfonate.
Notes: Drugs with a new additional indication for the treatment of systemic sclerosis associated interstitial lung disease. [Orphan drug]